PMDA approval: Tasigna Capsules 200 mg
Applicant: Novartis Pharma K.K. Approval date: 2009-01-21. Type: New approval. Active ingredient(s): Nilotinib hydrochloride hydrate.
Notes: A drug containing a new active ingredient indicated for the treatment of imatinib-resistant, chronic phase and accelerated phase chronic myelogenous leukemia. [Orphan drug]